PMDA approval: Opdivo Intravenous Infusion 20 mg Opdivo Intravenous Infusion 100 mg Opdivo Intravenous Infusion 120 mg Opdivo Intravenous Infusion 240 mg
Applicant: Ono Pharmaceutical Co., Ltd. Approval date: 2024-12-27. Type: Partial change. Active ingredient(s): Nivolumab (genetical recombination).
Notes: Drugs with a new indication and a new dosage for the treatment of unresectable urothelial carcinoma.